PMDA approval: INOflo for Inhalation 800 ppm
Applicant: INO Therapeutics LLC. Approval date: 2015-08-24. Type: Partial change. Active ingredient(s): Nitric oxide.
Notes: A drug with a new additional indication and a new dosage for the improvement of pulmonary hypertension in the perioperative period of cardiac surgery. [Orphan drug]